PMDA approval: Onepal No. 1 Injection Onepal No. 2 Injection
Applicant: AY Pharmaceuticals Co., Ltd. Approval date: 2018-03-23. Type: New approval. Active ingredient(s): N/A for this combination drug.
Notes: Combination prescription drugs with similar formulations indicated for the supplementation of water, electrolytes, amino acids, calories, vitamins, zinc, iron, copper, manganese, and iodine in patients for whom receiving oral or enteral nutrition is impossible or insufficient and the total parenteral nutrition is the only choice.